PMDA approval: Feron for Injection 6 MIU Feron for Injection 3 MIU Feron for Injection 1 MIU
Applicant: Toray Industries, Inc. Approval date: 2009-10-16. Type: Partial change. Active ingredient(s): Interferon beta.
Notes: Drugs with a new additional indication and a new dosage for the improvement of viraemia in chronic hepatitis C on concomitant administration with ribavirin. Rebetol Capsule 200 mg (Schering-Plough K.K.) Change Ribavirin A drug with a new additional indication and a new dosage for the improvement of viraemia in chronic hepatitis C on concomitant administration with interferon beta.